PMDA approval: Remitoro for Intravenous Drip Infusion 300 μg
Applicant: Eisai Co., Ltd. Approval date: 2021-03-23. Type: New approval. Active ingredient(s): Denileukin diftitox (genetical recombination).
Notes: A drug with a new active ingredient indicated for the treatment of relapsed or refractory peripheral T-cell lymphoma and relapsed or refractory cutaneous T-cell lymphoma.